PMDA approval: Givlaari Subcutaneous Injection 189 mg
Applicant: Alnylam Japan K.K. Approval date: 2021-06-23. Type: New approval. Active ingredient(s): Givosiran sodium.
Notes: A drug with a new active ingredient indicated for the treatment of acute hepatic porphyria. [Orphan drug]